PMDA approval: Nexium Capsules 10 mg Nexium Capsules 20 mg
Applicant: AstraZeneca K.K. Approval date: 2012-06-22. Type: Partial change. Active ingredient(s): Esomeprazole magnesium hydrate.
Notes: Drugs with a new additional indication and a new dosage for the prevention of recurrence of gastric ulcer or duodenal ulcer in patients treated with low-dose aspirin.